PMDA approval: Sargmalin for Inhalation 250 μg
Applicant: Nobelpharma Co., Ltd. Approval date: 2024-03-26. Type: New approval. Active ingredient(s): Sargramostim (genetical recombination).
Notes: A drug with a new active ingredient indicated for the treatment of autoimmune pulmonary alveolar proteinosis. [Orphan drug]